PMDA approval: Setaneo Ophthalmic Solution 0.002%
Applicant: Santen Pharmaceutical Co., Ltd. Approval date: 2025-08-25. Type: New approval. Active ingredient(s): Sepetaprost.
Notes: A drug with a new active ingredient indicated for the treatment of glaucoma and ocular hypertension.